PMDA approval: Verquvo Tablets 2.5 mg Verquvo Tablets 5 mg Verquvo Tablets 10 mg
Applicant: Bayer Yakuhin, Ltd. Approval date: 2021-06-23. Type: New approval. Active ingredient(s): Vericiguat.
Notes: Drugs with a new active ingredient indicated for the treatment of chronic heart failure (for use only in patients receiving standard treatment of chronic heart failure).